PMDA approval: Trastuzumab BS for I.V. Infusion 60 mg "NK" Trastuzumab BS for I.V. Infusion 150 mg "NK" (Nippon Kayaku Co., Ltd.) Trastuzumab BS for I.V. Infusion 60 mg "CTH" Trastuzumab BS for I.V. Infusion 150 mg "CTH"
Applicant: Celltrion Inc. Approval date: 2018-03-23. Type: New approval. Active ingredient(s): Trastuzumab (genetical recombination) [trastuzumab biosimilar 1].
Notes: Follow-on biologics indicated for the treatment of unresectable advanced or recurrent gastric cancer with HER2 overexpression.